PMDA approval: Opdivo Intravenous Infusion 20 mg Opdivo Intravenous Infusion 100 mg
Applicant: Ono Pharmaceutical Co., Ltd. Approval date: 2017-09-22. Type: Partial change. Active ingredient(s): Nivolumab (genetical recombination).
Notes: Drugs with a new additional indication for the treatment of unresectable advanced or recurrent gastric cancer which has progressed after cancer chemotherapy. [Priority review]